PMDA approval: Elplat I.V. Infusion Solution 50 mg Elplat I.V. Infusion Solution 100 mg Elplat for Injection 50 mg Elplat for Injection 100 mg
Applicant: Yakult Honsha Co., Ltd. Approval date: 2009-08-20. Type: New approval. Active ingredient(s): Oxaliplatin.
Notes: Drugs with a new additional indication and a new dosage in an additional dosage form (Elplat I.V. Infusion Solution 50 mg and Elplat I.V. Infusion Solution 100 mg) for post-operative adjuvant chemotherapy for colon cancer. [Priority review]